PMDA approval: Empliciti for I.V. Infusion 300 mg Empliciti for I.V. Infusion 400 mg
Applicant: Bristol-Myers Squibb Company. Approval date: 2016-09-28. Type: New approval. Active ingredient(s): Elotuzumab (genetical recombination).
Notes: Drugs with a new active ingredient indicated for the treatment of relapsed or refractory multiple myeloma. [Orphan drug]